PMDA approval: Ceprotin for Intravenous Injection 1000 IU
Applicant: Takeda Pharmaceutical Company Limited. Approval date: 2024-03-26. Type: New approval. Active ingredient(s): Human protein C.
Notes: A drug with a new active ingredient indicated for the treatment of venous thromboembolism and purpura fulminans and for the control of thrombophilia, in patients with congenital protein C deficiency.